PMDA approval: Spinraza Intrathecal injection 12 mg
Applicant: Biogen Japan Ltd. Approval date: 2017-07-03. Type: New approval. Active ingredient(s): Nusinersen sodium.
Notes: A drug with a new active ingredient indicated for the treatment of infantile spinal muscular atrophy. [Orphan drug]